PMDA approval: Xevudy for Intravenous Injection 500 mg
Applicant: GlaxoSmithKline K.K. Approval date: 2021-09-27. Type: New approval. Active ingredient(s): Sotrovimab (genetical recombination).
Notes: A drug with a new active ingredient indicated for the treatment of disease caused by SARS-CoV-2 infection (COVID-19). [Drug pertaining to Special Approval for Emergency]